PMDA approval: Linzess Tablets 0.25 mg
Applicant: Astellas Pharma Inc. Approval date: 2016-12-19. Type: New approval. Active ingredient(s): Linaclotide.
Notes: A drug with a new active ingredient indicated for the treatment of irritable bowel syndrome with constipation.